PMDA approval: Oxis 9 μg Turbuhaler 28 doses Oxis 9 μg Turbuhaler 60 doses
Applicant: AstraZeneca K.K. Approval date: 2012-06-29. Type: New approval. Active ingredient(s): Formoterol fumarate hydrate.
Notes: Drugs with a new indication and a new dosage for the alleviation of various symptoms due to airway obstructive impairment in chronic obstructive pulmonary diseases (chronic bronchitis and emphysema).